PMDA approval: Nasanyl Nasal Spray 0.2%
Applicant: Pfizer Japan Inc. Approval date: 2022-08-24. Type: Partial change. Active ingredient(s): Nafarelin acetate hydrate.
Notes: A drug with a new indication and a new dosage for the prevention of premature ovulation in assisted reproductive technology. [Public knowledge-based application after PAFSC's preliminary assessment]